PMDA approval: Erleada Tablets 60 mg
Applicant: Janssen Pharmaceutical K.K. Approval date: 2020-05-29. Type: Partial change. Active ingredient(s): Apalutamide.
Notes: A drug with a new indication for the treatment of metastatic prostate cancer.